PMDA approval: Dafclir Tablets 200 mg
Applicant: Astellas Pharma Inc. Approval date: 2018-07-02. Type: New approval. Active ingredient(s): Fidaxomicin.
Notes: A drug with a new active ingredient indicated for the treatment of infectious enteritis (including pseudomembranous colitis) caused by C. difficile.